PMDA approval: Lamictal Tablets 25 mg Lamictal Tablets 100 mg
Applicant: GlaxoSmithKline K.K. Approval date: 2011-07-01. Type: Partial change. Active ingredient(s): Lamotrigine.
Notes: Drugs with a new additional indication and a new dosage for the prevention of recurrence/relapse of mood episodes in patients with bipolar disorder.